PMDA approval: Ovisot for Injection 0.1 g
Applicant: Daiichi Sankyo Company, Limited. Approval date: 2017-08-25. Type: Partial change. Active ingredient(s): Acetylcholine chloride.
Notes: A drug with a new route of administration indicated for the induction of coronary spasm in a drug-induced coronary spasm provocation test during coronary angiography. [Public knowledge-based application after PAFSC's preliminary assessment]